PMDA approval: Maviret Combination Tablets
Applicant: AbbVie GK. Approval date: 2019-08-22. Type: Partial change. Active ingredient(s): Glecaprevir hydrate/Pibrentasvir.
Notes: A drug with a new additional pediatric dosage indicated for the improvement of viremia in patients with chronic hepatitis C or compensated cirrhosis type C. [Priority review]